PMDA approval: Sovaldi Tablets 400 mg
Applicant: Gilead Sciences, Inc. Approval date: 2015-03-26. Type: New approval. Active ingredient(s): Sofosbuvir.
Notes: A drug with a new active ingredient indicated for the improvement of viremia in patients with chronic hepatitis C or compensated cirrhosis type C in serogroup 2 (genotype 2). [Priority review]